PMDA approval: Genotropin 5.3 mg Genotropin MiniQuick S.C. Inj. 0.4 mg Genotropin MiniQuick S.C. Inj. 0.6 mg Genotropin MiniQuick S.C. Inj. 0.8 mg Genotropin MiniQuick S.C. Inj. 1.0 mg Genotropin MiniQuick S.C. Inj. 1.4 mg Genotropin Inj. 12 mg
Applicant: Pfizer Japan Inc. Approval date: 2008-10-16. Type: Partial change. Active ingredient(s): Somatropin (genetical recombination).
Notes: Drugs with a new indication and a new dosage for the treatment of dwarfism with no epiphyseal closure in patients born small-for-gestational age (SGA).